PMDA approval: Menactra Intramuscular Injection
Applicant: Sanofi K.K. Approval date: 2014-07-04. Type: New approval. Active ingredient(s): Meningococcal quadrivalent vaccine (diphtheria toxoid conjugate).
Notes: A drug with a new active ingredient indicated for the prophylaxis of invasive meningococcal disease. (serotypes A, C, Y, and W-135).